PMDA approval: Biktarvy Combination Tablets
Applicant: Gilead Sciences K.K. Approval date: 2019-03-26. Type: New approval. Active ingredient(s): Bictegravir sodium/ Emtricitabine/Tenofovir alafenamide fumarate.
Notes: A new combination drug with a new active ingredient indicated for the treatment of HIV-1 infection. [Orphan drug]